PMDA approval: Humalog Mix 50 Cart Humalog Mix 50 Kit Humalog Mix 50 MirioPen
Applicant: Eli Lilly Japan K.K. Approval date: 2009-08-20. Type: Partial change. Active ingredient(s): Insulin lispro (genetical recombination).
Notes: Drugs with a new dosage indicated for the treatment of diabetes mellitus where insulin therapy is indicated.